PMDA approval: Soliqua Injection Solostar
Applicant: Sanofi K.K. Approval date: 2020-03-25. Type: New approval. Active ingredient(s): Insulin glargine (genetical recombination)/ Lixisenatide.
Notes: A new combination drug indicated for the treatment of type 2 diabetes mellitus in cases where insulin therapy is indicated.